PMDA approval: Velcade Injection 3 mg
Applicant: Janssen Pharmaceutical K.K. Approval date: 2011-09-16. Type: Partial change. Active ingredient(s): Bortezomib.
Notes: A drug with a new indication and a new dosage for the treatment of multiple myeloma. [Orphan drug]